PMDA approval: Vabysmo Solution for Intravitreal Injection 120 mg/mL Vabysmo Kit for Intravitreal Injection 120 mg/mL
Applicant: Chugai Pharmaceutical Co., Ltd. Approval date: 2025-05-19. Type: Partial change. Active ingredient(s): Faricimab (genetical recombination).
Notes: Drugs with a new indication and a new dosage for the treatment of choroidal neovascularization associated with angioid streaks. [Orphan drug]